PMDA approval: Predonine Tablets 5 mg (Shionogi & Co., Ltd.) Prednisolone Tablets 1 mg (Asahi Kasei) Prednisolone Tablets 5 mg (Asahi Kasei) (Asahi Kasei Pharma Corporation) Prednisolone Tablets 5 mg "Takeda" Prednisolone Powder 1% "Takeda"
Applicant: Takeda Pharmaceutical Company Limited. Approval date: 2011-09-16. Type: Partial change. Active ingredient(s): Prednisolone.
Notes: Drugs with a new indication for the treatment of multiple myeloma. [Expedited review]